PMDA approval: Entyvio Pens for S.C. Injection 108 mg Entyvio Syringes for S.C. Injection 108 mg
Applicant: Takeda Pharmaceutical Company Limited. Approval date: 2023-09-25. Type: Partial change. Active ingredient(s): Vedolizumab (genetical recombination).
Notes: Drugs with a new indication for the maintenance therapy of moderate to severe active Crohn’s disease (for use only in patients who have not sufficiently responded to conventional treatments).